PMDA approval: l'estrogel 0.06%
Applicant: Fuji Pharma Co., Ltd. Approval date: 2022-03-11. Type: Partial change. Active ingredient(s): Estradiol.
Notes: A drug with a new indication and a new dosage for adjusting the start timing of controlled ovarian stimulation in assisted reproductive technology and hormone replacement cycle in frozen-thawed embryo transfer. [Public knowledge-based application]